PMDA approval: Adalimumab BS Subcutaneous Injection 20 mg Syringe 0.2 mL "MA" Adalimumab BS Subcutaneous Injection 40 mg Syringe 0.4 mL "MA" Adalimumab BS Subcutaneous Injection 80 mg Syringe 0.8 mL "MA" Adalimumab BS Subcutaneous Injection 40 mg Pen 0.4 mL "MA"
Applicant: Mochida Pharmaceutical Co., Ltd. Approval date: 2021-03-23. Type: New approval. Active ingredient(s): Adalimumab (genetical recombination) [adalimumab biosimilar 3].
Notes: Follow-on biologics indicated for the treatment of rheumatoid arthritis (including the prevention of structural joint damage), and the treatment of the following diseases in patients who have not responded sufficiently to conventional treatments; polyarticular-course juvenile idiopathic arthritis, plaque psoriasis, psoriatic arthritis, pustular psoriasis, ankylosing spondylitis and intestinal Behçet's disease; the remission induction therapy and maintenance therapy for moderate to severe active Crohn's disease, and treatment of moderate to severe ulcerative colitis.